PMDA approval: Berinert P I.V. Injection 500
Applicant: CSL Behring K.K. Approval date: 2017-03-24. Type: Partial change. Active ingredient(s): Lyophilized human C1-inactivator concentrate.
Notes: A drug with a new additional indication and a new dosage for inhibiting onset of acute attacks of hereditary angioedema caused by invasive procedures. [Public knowledge-based application after PAFSC's preliminary assessment]